PMDA approval: Lyfnua Tablets 45 mg
Applicant: MSD K.K. Approval date: 2022-01-20. Type: New approval. Active ingredient(s): Gefapixant citrate.
Notes: A drug with a new active ingredient indicated for the treatment of refractory chronic cough.